PMDA approval: Lenvima Capsule 4 mg Lenvima Capsule 10 mg
Applicant: Eisai Co., Ltd. Approval date: 2021-03-23. Type: Partial change. Active ingredient(s): Lenvatinib mesilate.
Notes: Drugs with a new indication for the treatment of unresectable thymic carcinoma. [Orphan drug]